PMDA approval: Concerta Tablets 18 mg Concerta Tablets 27 mg
Applicant: Janssen Pharmaceutical K.K. Approval date: 2013-12-20. Type: Partial change. Active ingredient(s): Methylphenidate hydrochloride.
Notes: Drugs with a new additional indication and a new dosage for the treatment of attention-deficit/hyperactivity disorder (AD/HD) in adults.